PMDA approval: ABILIFY Prolonged Release Aqueous Suspension for IM Injection 300 mg ABILIFY Prolonged Release Aqueous Suspension for IM Injection 400 mg ABILIFY Prolonged Release Aqueous Suspension for IM Injection 300 mg Syringes ABILIFY Prolonged Release Aqueous Suspension for IM Injection 400 mg Syringes
Applicant: Otsuka Pharmaceutical Co., Ltd. Approval date: 2020-09-25. Type: Partial change. Active ingredient(s): Aripiprazole hydrate.
Notes: Drugs with a new indication for the prevention of recurrence/relapse of mood episodes in patients with bipolar I disorder.